PMDA approval: Precedex Intravenous Solution 200 µg "Hospira" (Hospira Japan Co., Ltd.) Precedex Intravenous Solution 200 µg "Maruishi"
Applicant: Maruishi Pharmaceutical Co., Ltd. Approval date: 2013-06-14. Type: Partial change. Active ingredient(s): Dexmedetomidine hydrochloride.
Notes: Drugs with a new additional indication and a new dosage for sedation of non-intubated patients during surgical and other procedures under local anesthesia.